PMDA approval: Obizur Intravenous Injection 500
Applicant: Takeda Pharmaceutical Company Limited. Approval date: 2024-03-26. Type: New approval. Active ingredient(s): Susoctocog alfa (genetical recombination).
Notes: A drug with a new active ingredient indicated for the control of bleeding episodes in patients with acquired hemophilia A. [Orphan drug]